PMDA approval: YazFlex Combination Tablets
Applicant: Bayer Yakuhin Ltd. Approval date: 2016-12-19. Type: New approval. Active ingredient(s): Drospirenone /Ethinylestradiol betadex.
Notes: A drug with new indications and a new dosage in an additional dosage form indicated for the alleviation of pain associated with endometriosis, and dysmenorrhea.